PMDA approval: Adriacin Injection 10 Adriacin Injection 50
Applicant: Sandoz Pharma K.K. Approval date: 2022-11-24. Type: Partial change. Active ingredient(s): Doxorubicin hydrochloride.
Notes: Drugs with a new dosage indicated for the treatment of breast cancer (preoperative or postoperative chemotherapy in patients with operable breast cancer). [Public knowledge-based application]